PMDA approval: Rifxima Tablets 200 mg
Applicant: Aska Pharmaceutical Co., Ltd. Approval date: 2016-09-28. Type: New approval. Active ingredient(s): Rifaximin.
Notes: A drug with a new active ingredient indicated for the improvement of hyperammonemia in patients with hepatic encephalopathy. [Orphan drug]